PMDA approval: Aflibercept BS Solution for Intravitreal Injection 40 mg/mL "GRP"
Applicant: Global Regulatory Partners GK. Approval date: 2024-06-24. Type: New approval. Active ingredient(s): Aflibercept (genetical recombination) [aflibercept biosimilar 1].
Notes: A biosimilar indicated for the treatment of macular edema following retinal vein occlusion, choroidal neovascularization in patients with pathologic myopia, and diabetic macular edema.